PMDA approval: Gracevit Tablets 50 mg Gracevit Fine Granules 10%
Applicant: Daiichi Sankyo Company, Limited. Approval date: 2011-08-17. Type: Partial change. Active ingredient(s): Sitafloxacin hydrate.
Notes: Drugs with a new dosage indicated for the treatment of pharyngitis and laryngitis, tonsillitis (including peritonsillitis and peritonsillar abscess), acute bronchitis, pneumonia, secondary infection of chronic respiratory disease, cystitis, pyelonephritis, urethritis, cervicitis, otitis media, sinusitis, periodontal inflammation, pericoronitis, and jaw inflammation.